PMDA approval: Elplat for Injection 50 mg Elplat for Injection 100 mg Elplat I.V. Infusion Solution 50 mg Elplat I.V. Infusion Solution 100 mg
Applicant: Yakult Honsha Co., Ltd. Approval date: 2011-11-25. Type: Partial change. Active ingredient(s): Oxaliplatin.
Notes: Drugs with a new dosage for post-operative adjuvant chemotherapy for colon cancer.